PMDA approval: Vonvendi Intravenous 1300
Applicant: Shire Japan K.K. Approval date: 2020-03-25. Type: New approval. Active ingredient(s): Vonicog alfa (genetical recombination).
Notes: A drug with a new active ingredient indicated for the control of bleeding tendency in patients with von Willebrand disease (VWD). [Orphan drug]